PMDA approval: (1) Koselugo Granules 5 mg (2) Koselugo Granules 7.5 mg (3) Koselugo Capsules 10 mg (4) Koselugo Capsules 25 mg
Applicant: Alexion Pharma GK. Approval date: 2025-09-19. Type: New approval. Active ingredient(s): Selmetinib sulfate (1) (2).
Notes: Drugs with a new dosage in an additional dosage form indicated for the treatment of plexiform neurofibroma in patients with neurofibromatosis type 1. (3) (4) Drugs with a new dosage indicated for the treatment of plexiform neurofibroma in patients with neurofibromatosis type 1. [Orphan drug]